PMDA approval: Sutent Capsule 12.5 mg
Applicant: Pfizer Japan Inc. Approval date: 2012-08-10. Type: Partial change. Active ingredient(s): Sunitinib malate.
Notes: A drug with a new additional indication and a new dosage for pancreatic neuroendocrine tumour. [Orphan drug]